PMDA approval: Zokinvy Capsules 50 mg Zokinvy Capsules 75 mg
Applicant: AnGes, Inc. Approval date: 2024-01-18. Type: New approval. Active ingredient(s): Lonafarnib.
Notes: Drugs with a new active ingredient indicated for the treatment of Hutchinson-Gilford Progeria syndrome and processing-deficient progeroid laminopathies. [Orphan drug]